PMDA approval: Vfend Tablets 50 mg Vfend Tablets 200 mg Vfend for Intravenous Use 200 mg Vfend Dry Syrup 2800 mg
Applicant: Pfizer Japan Inc. Approval date: 2014-09-26. Type: Partial change. Active ingredient(s): Voriconazole.
Notes: Drugs with new additional pediatric dosages, and a drug with a new additional pediatric dosage in an additional dosage form of dry syrup indicated for the treatment of severe or refractory fungus infection.